PMDA approval: Treprost Inhalation Solution 1.74 mg
Applicant: Mochida Pharmaceutical Co., Ltd. Approval date: 2024-09-24. Type: Partial change. Active ingredient(s): Treprostinil.
Notes: A drug with a new indication and a new dosage for the treatment of pulmonary hypertension associated with interstitial lung disease. [Orphan drug]